PMDA approval: Diflucan Intravenous Solution 50 mg Diflucan Intravenous Solution 100 mg Diflucan Intravenous Solution 200 mg Diflucan Capsules 50 mg Diflucan Capsules 100 mg
Applicant: Pfizer Japan Inc. Approval date: 2011-11-25. Type: Partial change. Active ingredient(s): Fluconazole.
Notes: Drugs with a new additional pediatric dosage and a new additional indication for the prevention of deep mycosis in patients with hematopoietic stem cell transplantation. [Public knowledge-based application after PAFSC's preliminary assessment]